PMDA approval: Marizev Tablets 25 mg Marizev Tablets 12.5 mg
Applicant: MSD K.K. Approval date: 2015-09-28. Type: New approval. Active ingredient(s): Omarigliptin.
Notes: Drugs with a new active ingredient indicated for the treatment of type 2 diabetes mellitus.